PMDA approval: Minnebro Tablets 1.25 mg Minnebro Tablets 2.5 mg Minnebro Tablets 5 mg
Applicant: Daiichi Sankyo Company, Limited. Approval date: 2019-01-08. Type: New approval. Active ingredient(s): Esaxerenone.
Notes: Drugs with a new active ingredient indicated for the treatment of hypertension.